PMDA approval: Growject S.C. Injection 6 mg Growject S.C. Injection 12 mg
Applicant: JCR Pharmaceuticals Co., Ltd. Approval date: 2023-06-26. Type: Partial change. Active ingredient(s): Somatropin (genetical recombination).
Notes: Drugs with a new indication for the treatment of short stature in SHOX deficiency prior to epiphyseal closure.